PMDA approval: Tresiba Flex Touch Tresiba Penfill
Applicant: Novo Nordisk Pharma Ltd. Approval date: 2012-09-28. Type: New approval. Active ingredient(s): Insulin degludec (genetical recombination).
Notes: Drugs with a new active ingredient indicated for the treatment of diabetes mellitus in cases where insulin therapy is indicated.